PMDA approval: Hiyasta Tablets 10 mg
Applicant: Huya Japan G.K. Approval date: 2021-11-25. Type: Partial change. Active ingredient(s): Tucidinostat.
Notes: A drug with a new indication for the treatment of relapsed or refractory peripheral T-cell lymphoma. [Orphan drug]